PMDA approval: Welireg Tablets 40 mg
Applicant: MSD K.K. Approval date: 2025-06-24. Type: New approval. Active ingredient(s): Belzutifan.
Notes: (1) A drug with a new active ingredient indicated for the treatment of unresectable or metastatic renal cell carcinoma that has progressed after cancer chemotherapy. (2) A drug with a new active ingredient indicated for the treatment of von Hippel-Lindau disease-associated tumors. [Orphan Drug (only for [2])]